PMDA approval: Norvasc Tablets 2.5 mg Norvasc Tablets 5 mg Norvasc OD Tablets 2.5 mg Norvasc OD Tablets 5 mg (Pfizer Japan Inc.) Amlodin Tablets 2.5 mg Amlodin Tablets 5 mg Amlodin OD Tablets 2.5 mg Amlodin OD Tablets 5 mg
Applicant: Dainippon Sumitomo Pharma Co., Ltd. Approval date: 2009-02-23. Type: Partial change. Active ingredient(s): Amlodipine besilate.
Notes: Drugs with a new dosage indicated for the treatment of hypertension and angina pectoris.